PMDA approval: Signifor LAR Kit for i.m. injection 10 mg Signifor LAR Kit for i.m. injection 30 mg Signifor LAR Kit for i.m. injection 20 mg Signifor LAR Kit for i.m. injection 40 mg
Applicant: Novartis Pharma K.K. Approval date: 2018-03-23. Type: New approval. Active ingredient(s): Pasireotide pamoate.
Notes: Drugs with a new additional indication and a new dosage and in additional dosage forms indicated for the treatment of Cushing’s disease (when surgical therapies are not sufficiently effective or are difficult to perform). [Orphan drug]